PMDA approval: Erbitux Injection 100 mg
Applicant: Merck KGaA. Approval date: 2008-07-16. Type: New approval. Active ingredient(s): Cetuximab (genetical recombination).
Notes: A drug containing a new active ingredient indicated for the treatment of EGFR-expressing, unresectable and advanced/recurrent colorectal carcinomas. [Priority review]